PMDA approval: Elrexfio S.C. Injection 44 mg Elrexfio S.C. Injection 76 mg
Applicant: Pfizer Japan Inc. Approval date: 2024-03-26. Type: New approval. Active ingredient(s): Elranatamab (genetical recombination).
Notes: Drugs with a new active ingredient indicated for the treatment of relapsed or refractory multiple myeloma (for use only if refractory or intolerant to standard therapies). [Orphan drug]